PMDA approval: Julina Tablets 0.5 mg
Applicant: Bayer Yakuhin, Ltd. Approval date: 2022-03-11. Type: Partial change. Active ingredient(s): Estradiol.
Notes: A drug with a new indication and a new dosage for adjusting the start timing of controlled ovarian stimulation in assisted reproductive technology and hormone replacement cycle in frozen-thawed embryo transfer. [Public knowledge-based application]